PMDA approval: Vidaza for Injection 100 mg
Applicant: Nippon Shinyaku Co., Ltd. Approval date: 2021-03-23. Type: Partial change. Active ingredient(s): Azacitidine.
Notes: A drug with a new indication for the treatment of acute myeloid leukemia. [Expedited review]